PMDA approval: Fludara for IV Inj.50 mg
Applicant: Bayer Yakuhin, Ltd. Approval date: 2008-05-20. Type: Partial change. Active ingredient(s): Fludarabine phosphate.
Notes: A drug with a new indication and a new dosage for use as a conditioning prior to allogeneic hematopoietic stem cell transplantation in the following diseases: acute myeloid leukemia, myelodysplastic syndrome, chronic myeloid leukemia, chronic lymphocytic leukemia, malignant lymphoma, and multiple myeloma.